PMDA approval: NovoSeven for Injection 1.2 mg NovoSeven for Injection 4.8 mg NovoSeven HI for Intravenous Injection 1 mg NovoSeven HI for Intravenous Injection 2 mg NovoSeven HI for Intravenous Injection 5 mg
Applicant: Novo Nordisk Pharma Ltd. Approval date: 2011-06-16. Type: Partial change. Active ingredient(s): Eptacog alfa (activated) (genetical recombination).
Notes: Drugs with a new additional indication and a new dosage for inhibition of bleeding tendency in patients with Glanzmann thrombasthenia with alloantibodies against platelet, and with past or present refractoriness to platelet transfusions. [Public knowledge-based application after PAFSC's preliminary assessment]